PMDA approval: Diagnogreen for Injection 25 mg
Applicant: Daiichi Sankyo Company, Limited. Approval date: 2018-07-02. Type: Partial change. Active ingredient(s): Indocyanine green.
Notes: A drug with a new additional indication and a new dosage for the evaluation of vascular and tissue blood flow. [Public knowledge-based application after PAFSC's preliminary assessment]